PMDA approval: Lasvic Tablets 75 mg
Applicant: Kyorin Pharmaceutical Co., Ltd. Approval date: 2019-09-20. Type: New approval. Active ingredient(s): Lascufloxacin hydrochloride.
Notes: A drug with a new active ingredient indicated for the treatment of laryngopharyngitis, tonsillitis (including peritonsillitis and peritonsillar abscess), acute bronchitis, pneumonia, secondary infection of chronic respiratory disease, otitis media, and sinusitis caused by Lasvic-sensitive Staphylococcus spp., Streptococcus spp., Streptococcus pneumoniae, Moraxella (Branhamella) catarrhalis, Klebsiella spp., Enterobacter spp., Haemophilus influenzae, Legionella pneumophila, Prevotella spp., and Mycoplasma pneumoniae.